PMDA approval: Micamlo Combination Tablets BP
Applicant: Nippon Boehringer Ingelheim Co., Ltd. Approval date: 2012-12-21. Type: New approval. Active ingredient(s): Telmisartan/amlodipine besilate.
Notes: A drug with a new dosage in an additional dosage form for the treatment of hypertension.